PMDA approval: Keytruda Injection 100 mg Keytruda Injection 20 mg
Applicant: MSD K.K. Approval date: 2018-12-21. Type: Partial change. Active ingredient(s): Pembrolizumab (genetical recombination).
Notes: Drugs with new indications and a new dosage for the treatment of: (1) advanced or recurrent microsatellite instability-high (MSI-High) solid tumors that have progressed after cancer chemotherapy (for use only if refractory or intolerant to standard therapies), (2) melanoma, and (3) unresectable advanced or recurrent non-small cell lung cancer. [(1) Conditional early approval, (2) Orphan drug, (3) Priority review]